PMDA approval: FP-OD Tablet 2.5
Applicant: FP Pharmaceutical Corporation. Approval date: 2015-12-21. Type: Partial change. Active ingredient(s): Selegiline hydrochloride.
Notes: A drug with a revised indication and a new dosage for the treatment of Parkinson's disease (When used in combination with levodopa-containing products; patients are classified as Stage I to IV on the Hoehn and Yahr scale, and when not used in combination with levodopa-containing products; patients are classified as Stage I to III on the Hoehn and Yahr scale).